PMDA approval: Jakavi Tablets 5 mg
Applicant: Novartis Pharma K.K. Approval date: 2015-09-24. Type: Partial change. Active ingredient(s): Ruxolitinib phosphate.
Notes: A drug with a new additional indication and a new dosage for the treatment of polycythemia vera (use only when conventional therapies are not sufficiently effective or inappropriate).